PMDA approval: Ultomiris for Intravenous Infusion 300 mg Ultomiris for Intravenous Infusion 300 mg/3 mL Ultomiris for Intravenous Infusion 1100 mg/11 mL
Applicant: Alexion Pharma GK. Approval date: 2022-08-24. Type: Partial change. Active ingredient(s): Ravulizumab (genetical recombination).
Notes: Drugs with a new indication for the treatment of generalized myasthenia gravis (for use only in patients whose symptoms cannot be sufficiently controlled with high-dose intravenous immunoglobulin therapy or plasmapheresis).